PMDA approval: Relvar 100 Ellipta 14 dose Relvar 100 Ellipta 30 dose
Applicant: GlaxoSmithKline K.K. Approval date: 2016-12-02. Type: Partial change. Active ingredient(s): Vilanterol trifenatate/ Fluticasone furoate.
Notes: Drugs with a new additional indication and a new dosage for the relief of symptoms in patients with chronic obstructive pulmonary disease (chronic bronchitis, emphysema) (who require a combination therapy with an inhaled corticosteroid and a long-acting beta-2 agonist).